PMDA approval: Maviret Combination Tablets Maviret Combination Granules for Pediatric Use
Applicant: AbbVie GK. Approval date: 2022-06-20. Type: Partial change. Active ingredient(s): Glecaprevir hydrate/Pibrentasvir.
Notes: A drug with an additional pediatric dosage (aged 3 years to 11 years) and a drug with an additional pediatric dosage (aged 3 years to 11 years) in an additional dosage form, indicated for the improvement of viremia in patients with chronic hepatitis C or compensated cirrhosis type C.